PMDA approval: Rasimlo Combination Tablet LD Rasimlo Combination Tablet HD
Applicant: Novartis Pharma K.K. Approval date: 2014-03-24. Type: New approval. Active ingredient(s): Aliskiren fumarate/amlodipine besylate.
Notes: New combination drugs indicated for the treatment of hypertension.